PMDA approval: Adoair 100 Diskus Adoair 250 Diskus Adoair 500 Diskus
Applicant: GlaxoSmithKline K.K. Approval date: 2007-04-18. Type: New approval. Active ingredient(s): Salmeterol xinafoate/Fluticasone propionate.
Notes: A new combination drug indicated for the treatment of asthma (in the case where a combination of inhaled corticosteroids and inhaled long-acting β2 agonists is necessary).